PMDA approval: Abstral Sublingual Tablet 100 μg Abstral Sublingual Tablet 200 μg Abstral Sublingual Tablet 400 μg
Applicant: Kyowa Hakko Kirin Co., Ltd. Approval date: 2013-09-20. Type: New approval. Active ingredient(s): Fentanyl citrate.
Notes: Drugs with a new dosage in a new dosage form indicated for analgesia of breakthrough pain in patients with cancer receiving a potent opioid analgesic at fixed time.